PMDA approval: Emulsion-adjuvanted Cell-culture Derived Influenza HA Vaccine (prototype) for Intramuscular Injection “Kaketsuken”
Applicant: Kaketsuken [The Chemo-Sero-Therapeutic Research Institute]. Approval date: 2018-03-23. Type: Partial change. Active ingredient(s): Emulsion-adjuvanted cell-culture derived influenza HA vaccine (prototype).
Notes: A drug with a new additional pediatric dosage indicated for the prevention of pandemic influenza. [Orphan drug]